PMDA approval: Thyradin-S Intravenous Injection 200 μg
Applicant: Aska Pharmaceutical Co., Ltd. Approval date: 2020-01-23. Type: New approval. Active ingredient(s): Levothyroxine sodium hydrate.
Notes: A drug with a new route of administration indicated for the treatment of myxedema coma and hypothyroidism (for hypothyroidism, only in patients ineligible for oral levothyroxine sodium therapy).